PMDA approval: Irribow Tablets 2.5 μg Irribow Tablets 5 μg Irribow OD Tablets 2.5 μg Irribow OD Tablets 5 μg
Applicant: Astellas Pharma Inc. Approval date: 2015-05-26. Type: Partial change. Active ingredient(s): Ramosetron hydrochloride.
Notes: Drugs with a new additional indication and a new dosage for the treatment of diarrhea-predominant irritable bowel syndrome in women.